PMDA approval: Rituxan Injection 10 mg/mL
Applicant: Zenyaku Kogyo Co., Ltd. Approval date: 2013-06-14. Type: Partial change. Active ingredient(s): Rituximab (genetical recombination).
Notes: A drug with new additional indications and a new dosage for the treatment of (1) CD 20-positive B-cell lymphoproliferative disorders in immunocompromised patients and (2) Wegener's granulomatosis and microscopic polyangiitis. [Public knowledge-based application after PAFSC's preliminary assessment]